PMDA approval: Leunase Injection 5000 Leunase Injection 10000
Applicant: Kyowa Hakko Kirin Co., Ltd. Approval date: 2013-02-21. Type: Partial change. Active ingredient(s): L-Asparaginase.
Notes: Drugs with a new route of administration. These drugs are indicated for acute leukemia (including blast crisis of chronic leukemia) and malignant lymphoma. [Public knowledge-based application after PAFSC's preliminary assessment]